PMDA approval: Adsorbed Pandemic Influenza Vaccine (H5N1) "Kaketsuken"
Applicant: Kaketsuken [The Chemo-Sero-Therapeutic Research Institute]. Approval date: 2010-10-27. Type: New approval. Active ingredient(s): Inactivated influenza virus, H5N1 (whole virion).
Notes: A drug with a new active ingredient indicated for the prevention of pandemic influenza (H5N1). [Orphan drug]